PMDA approval: (1) Talzenna Capsules 0.1 mg (2) Talzenna Capsules 0.25 mg (3) Talzenna Capsules 1 mg
Applicant: Pfizer Japan Inc. Approval date: 2024-01-18. Type: New approval. Active ingredient(s): Talazoparib tosilate.
Notes: (1) A drug with a new active ingredient indicated for the treatment of BRCA mutation-positive, metastatic castration-resistant prostate cancer. (2) A drug with a new active ingredient indicated for the treatment of BRCA mutation-positive, metastatic castration-resistant prostate cancer and unresectable or recurrent BRCA mutation-positive and HER2-negative breast cancer in patients who have previously been treated with chemotherapy. (3) A drug with a new active ingredient indicated for the treatment of unresectable or recurrent BRCA mutation-positive and HER2-negative breast cancer in patients who have previously been treated with chemotherapy.